PMDA approval: Iclusig Tablets 15 mg
Applicant: Otsuka Pharmaceutical Co., Ltd. Approval date: 2016-09-28. Type: New approval. Active ingredient(s): Ponatinib hydrochloride.
Notes: A drug with a new active ingredient indicated for the treatment of chronic myelogenous leukemia with resistance or intolerance to prior drug therapies, and recurrent or refractory Philadelphia chromosome-positive acute lymphoblastic leukemia. [Orphan drug]